PMDA approval: Remicade for I.V. Infusion 100
Applicant: Mitsubishi Tanabe Pharma Corporation. Approval date: 2009-07-07. Type: Partial change. Active ingredient(s): Infliximab (genetical recombination).
Notes: A drug with a new indication and a new dosage for the treatment of rheumatoid arthritis (including prevention of structural joint damage) in patients who have not sufficiently responded to conventional treatments. [Expedited review]